PMDA approval: Mulpleta Tablets 3 mg
Applicant: Shionogi & Co., Ltd. Approval date: 2015-09-28. Type: New approval. Active ingredient(s): Lusutrombopag.
Notes: A drug with a new active ingredient indicated for the improvement of thrombocytopenia in patients with chronic liver disease for whom an elective invasive procedure is planned.